PMDA approval: Penles Tape 18 mg
Applicant: Nitto Denko Corporation. Approval date: 2012-06-22. Type: Partial change. Active ingredient(s): Lidocaine.
Notes: A drug with a new additional indication and a new dosage for pain relief at the resection of molluscum contagiosum.